PMDA approval: 1) Wellnara Combination Tablets (Bayer Yakuhin, Ltd.) 2) Julina 0.5 mg
Applicant: Bayer Yakuhin, Ltd. Approval date: 2008-10-16. Type: New approval. Active ingredient(s): 1) Estradiol/levonorgestrel 2) Estradiol.
Notes: 1) A new combination drug and 2) a drug with a new indication and a new dosage for the treatment of postmenopausal osteoporosis.